PMDA approval: Patanol EX Ophthalmic Solution 0.2%
Applicant: Alcon Japan Ltd. Approval date: 2011-08-17. Type: New approval. Active ingredient(s): Olopatadine hydrochloride.
Notes: A drug with a new dosage and an additional dosage form indicated for the treatment of allergic conjunctivitis.